PMDA approval: Takelda Combination Tablets
Applicant: Takeda Pharmaceutical Company Limited. Approval date: 2014-03-24. Type: New approval. Active ingredient(s): Lansoprazole/aspirin.
Notes: A new combination drug indicated for the postoperative inhibition of thrombus and thrombus formation (limited to patients with previous history of gastric or duodenal ulcer) in patients with angina (chronic stable angina, unstable angina), myocardial infraction, ischemic cerebrovascular disorder (transient ischemic attack [TIA], cerebral infarction), and patients who have undergone coronary artery bypass grafting (CABG) or percutaneous transluminal coronary angioplasty (PTCA).